PMDA approval: Oncaspar I.V. Infusion 3750
Applicant: Nihon Servier Co., Ltd. Approval date: 2023-06-26. Type: New approval. Active ingredient(s): Pegaspargase.
Notes: A drug with a new active ingredient indicated for the treatment of acute lymphocytic leukemia and malignant lymphoma.